PMDA approval: Kenketu Glovenin-I-Nichiyaku
Applicant: Nihon Pharmaceutical Co., Ltd. Approval date: 2008-10-16. Type: Partial change. Active ingredient(s): Freeze-dried polyethylene glycol treated human normal immunoglobulin.
Notes: A drug with a new indication and a new dosage for the treatment of pemphigus (for use when steroid drugs are not sufficiently effective).